PMDA approval: Prograf Capsules 0.5 mg Prograf Capsules 1 mg Prograf Capsules 5 mg
Applicant: Astellas Pharma Inc. Approval date: 2009-07-07. Type: Partial change. Active ingredient(s): Tacrolimus hydrate.
Notes: Drugs with a new additional indication and a new dosage for the treatment of refractory (steroid-resistant/steroid-dependent) active ulcerative colitis (limited to moderate-to-severe cases).